PMDA approval: Tykerb Tablets 250 mg
Applicant: GlaxoSmithKline K.K. Approval date: 2015-11-20. Type: Partial change. Active ingredient(s): Lapatinib tosilate hydrate.
Notes: A drug with new dosages indicated for the treatment of inoperable or recurrent breast cancer with HER2 overexpression.